PMDA approval: Perjeta Intravenous Infusion 420 mg/14 mL
Applicant: Chugai Pharmaceutical Co., Ltd. Approval date: 2022-03-28. Type: Partial change. Active ingredient(s): Pertuzumab (genetical recombination).
Notes: A drug with a new indication and a new dosage for the treatment of unresectable advanced or recurrent HER2-positive colon or rectal cancer that has progressed after cancer chemotherapy.